PMDA approval: Mirena 52 mg
Applicant: Bayer Yakuhin, Ltd. Approval date: 2014-11-18. Type: Partial change. Active ingredient(s): Levonorgestrel.
Notes: A drug with a new additional indication for the treatment of dysmenorrhoea. [Public knowledge-based application after PAFSC's preliminary assessment]